PMDA approval: NovoRapid 100 U/ml
Applicant: Novo Nordisk Pharma Ltd. Approval date: 2012-06-22. Type: Partial change. Active ingredient(s): Insulin aspart (genetical recombination).
Notes: A drug with a new route of administration and a new dosage. This drug is indicated for the treatment of diabetes mellitus where insulin therapy is indicated.